PMDA approval: Etanercept BS 10 mg Syringe 1.0 mL for S.C. Inj. “TY” Etanercept BS 25 mg Syringe 0.5 mL for S.C. Inj. “TY” Etanercept BS 50 mg Syringe 1.0 mL for S.C. Inj. “TY” Etanercept BS 50 mg Pen 1.0 mL for S.C. Inj. “TY” (YL Biologics Limited) Etanercept BS 10 mg Syringe 1.0 mL for S.C. Inj. “Nichiiko” Etanercept BS 25 mg Syringe 0.5 mL for S.C. Inj. “Nichiiko” Etanercept BS 50 mg Syringe 1.0 mL for S.C. Inj. “Nichiiko” Etanercept BS 50 mg Pen 1.0 mL for S.C. Inj. “Nichiiko”
Applicant: Kyowa Pharmaceutical Industry Co., Ltd. Approval date: 2019-03-26. Type: New approval. Active ingredient(s): Etanercept (genetical recombination) [etanercept biosimilar 2].
Notes: Follow-on biologics indicated for the treatment of rheumatoid arthritis (including the prevention of structural joint damage) and polyarticular-course juvenile idiopathic arthritis in patients who have not responded sufficiently to conventional therapies.